PMDA approval: Myozyme 50 mg for Intravenous Infusion
Applicant: Genzyme Japan K.K. Approval date: 2007-04-18. Type: New approval. Active ingredient(s): Alglucosidase alfa (genetical recombination).
Notes: A drug containing a new active ingredient indicated for the treatment of type 2 glycogenosis. [Orphan drug]